PMDA approval: Imunomax-γ for Injection 50 Imunomax-γ for Injection 100
Applicant: Shionogi & Co., Ltd. Approval date: 2014-05-23. Type: Partial change. Active ingredient(s): Interferon gamma-1a (genetical recombination).
Notes: Drugs with new additional indications and a new dosage for the treatment of mycosis fungoides and Sézary syndrome. [Orphan drug]